PMDA approval: Hycamtin for Injection 1.1 mg
Applicant: Nippon Kayaku Co., Ltd. Approval date: 2011-02-23. Type: Partial change. Active ingredient(s): Nogitecan hydrochloride.
Notes: A drug with a new additional indication and a new dosage for the treatment of ovarian cancer which has progressed after cancer chemotherapy. [Public knowledge-based application after PAFSC's preliminary assessment]